PMDA approval: Tambocor Tablets 50 mg Tambocor Tablets 100 mg
Applicant: Eisai Co., Ltd. Approval date: 2010-05-13. Type: Partial change. Active ingredient(s): Flecainide acetate.
Notes: Drugs with a new additional indication and a new dosage for the treatment of tachyarrhythmia (paroxysmal atrial fibrillation/flutter, paroxysmal supraventricular tachycardia, ventricular tachycardia) in children. [Expedited review]